PMDA approval: Equa Tablets 50 mg
Applicant: Novartis Pharma K.K. Approval date: 2013-02-28. Type: Partial change. Active ingredient(s): Vildagliptin.
Notes: A drug with a new indication for the treatment of type 2 diabetes mellitus.